PMDA approval: [1] Neupro Patch 2.25 mg [2] Neupro Patch 4.5 mg [3] Neupro Patch 9 mg [4] Neupro Patch 13.5 mg
Applicant: Otsuka Pharmaceutical Co., Ltd. Approval date: 2012-12-25. Type: New approval. Active ingredient(s): Rotigotine [1] [2].
Notes: Drugs with a new active ingredient indicated for the treatment of Parkinson's disease and moderate to severe idiopathic restless legs syndrome. [3] [4] Drugs with a new active ingredient indicated for the treatment of Parkinson's disease.